PMDA approval: Colchicine Tablets 0.5 mg "Takata"
Applicant: Takata Pharmaceutical Co.,Ltd. Approval date: 2016-09-28. Type: Partial change. Active ingredient(s): Colchicine.
Notes: A drug with a new additional indication and a new dosage for the treatment of familial Mediterranean fever. [Public knowledge-based application after PAFSC's preliminary assessment]